PMDA approval: Takepron Capsules 15 Takepron Capsules 30 Takepron OD Tablets 15 Takepron OD Tablets 30 (Takeda Pharmaceutical Co., Ltd.) Omepral Tablets 10 mg Omepral Tablets 20 mg (AstraZeneca K.K.) Omeprazon Tablets 10 mg Omeprazon Tablets 20 mg (Mitsubishi Pharma Corporation) Pariet Tablets 10 mg (Eisai Co., Ltd.) Pasetocin Capsules Pasetocin Tablets 250 (Kyowa Hakko Kogyo Co., Ltd.) Sawacillin Capsules Sawacillin Tablets 250 (Takeda Pharmaceutical Co., Ltd.) Amolin Capsules 125 Amolin Capsules 250 10% Amolin Fine Granules (Astellas Pharma Inc.) Amopenixin Capsules 250 (Nipro Pharma Corporation) Flagyl (Shionogi & Co., Ltd.) Asuzol tablets
Applicant: Fuji Pharma Co., Ltd. Approval date: 2007-08-23. Type: Partial change. Active ingredient(s): Lansoprazole Omeprazole Rabeprazole sodium Amoxicillin Metronidazole.
Notes: Drugs with a new indication and dosage for use as a secondary eradication therapy for infection with Helicobacter pylori (3-drug combination therapy consisting of one-week dosing of a proton pump inhibitor (PPI), amoxicillin (AMPC), and metronidazole to patients in whom primary eradication therapy with PPI, AMPC, and clarithromycin was unsuccessful).